PMDA approval: [1] Predonine 10 mg [2] Predonine 20 mg [3] Predonine 50 mg [4] Predonine Tablets 5 mg (Shionogi & Co., Ltd.) [5] Prednisolone Tablets 1 mg (Asahi Kasei) [6] Prednisolone Tablets 5 mg (Asahi Kasei)
Applicant: Asahi Kasei Pharma Corporation. Approval date: 2013-09-13. Type: Partial change. Active ingredient(s): [1] - [3] Prednisolone sodium succinate [4] - [6] Prednisolone.
Notes: Drugs with a new additional indication and a new dosage for the treatment of acute-phase Kawasaki's disease (cases where the disease is severe and at risk for coronary artery disorder) ([1] - [6]) and Duchenne muscular dystrophy ([4] - [6]). Duchenne muscular dystrophy: [Public knowledge-based application after PAFSC's preliminary assessment]